PMDA approval: Onglyza Tablets 2.5 mg Onglyza Tablets 5 mg
Applicant: Otsuka Pharmaceutical Co., Ltd. Approval date: 2013-03-25. Type: New approval. Active ingredient(s): Saxagliptin hydrate.
Notes: Drugs with a new active ingredient indicated for the treatment of type 2 diabetes mellitus.